PMDA approval: Trastuzumab BS for Intravenous Drip Infusions 60 mg "Daiichi Sankyo" Trastuzumab BS for Intravenous Drip Infusions 150 mg "Daiichi Sankyo"
Applicant: Daiichi Sankyo Company, Limited. Approval date: 2018-09-21. Type: New approval. Active ingredient(s): Trastuzumab (genetical recombination) [trastuzumab biosimilar 2].
Notes: Follow-on biologics indicated for the treatment of breast cancer overexpressing HER2 and unresectable advanced or recurrent gastric cancer overexpressing HER2.